PMDA approval: Recomodulin Injection 12800
Applicant: Asahi Kasei Pharma Corporation. Approval date: 2008-01-25. Type: New approval. Active ingredient(s): Thrombomodulin alfa (genetical recombination).
Notes: A drug containing a new active ingredient indicated for the treatment of disseminated intravascular coagulation (DIC).